PMDA approval: Busulfex Injection 60 mg
Applicant: Otsuka Pharmaceutical Co., Ltd. Approval date: 2021-08-25. Type: Partial change. Active ingredient(s): Busulfan.
Notes: A drug with a new dosage indicated for the conditioning treatments prior to allogeneic hematopoietic stem cell transplantation, and autologous hematopoietic stem-cell transplantation for Ewing's sarcoma family of tumors and neuroblastoma. [Public knowledge-based application after PAFSC's preliminary assessment]